PMDA approval: Volibris Tablets 2.5 mg
Applicant: GlaxoSmithKline K.K. Approval date: 2010-07-23. Type: New approval. Active ingredient(s): Ambrisentan.
Notes: A drug with a new active ingredient indicated for the treatment of pulmonary arterial hypertension. [Orphan drug]